PMDA approval: NovoSeven HI Syringe for I.V. Injection 1 mg NovoSeven HI Syringe for I.V. Injection 2 mg NovoSeven HI Syringe for I.V. Injection 5 mg
Applicant: Novo Nordisk Pharma Ltd. Approval date: 2024-09-24. Type: Partial change. Active ingredient(s): Eptacog alfa (activated) (genetical recombination).
Notes: Drugs with a new indication and a new dosage for the control of bleeding tendency in patients with Glantzmann thrombasthenia. [Public knowledge-based application]